PMDA approval: Lorbrena Tablets 25 mg Lorbrena Tablets 100 mg
Applicant: Pfizer Japan Inc. Approval date: 2021-11-25. Type: Partial change. Active ingredient(s): Lorlatinib.
Notes: Drugs with a new indication for the treatment of unresectable or advanced/recurrent anaplastic lymphoma kinase (ALK) fusion gene-positive non-small cell lung cancer.